PMDA approval: Afinitor Tablets 5 mg
Applicant: Novartis Pharma K.K. Approval date: 2010-01-20. Type: New approval. Active ingredient(s): Everolimus.
Notes: A drug with a new additional indication and a new dosage for the treatment of unresectable or metastatic renal cell carcinoma. [Priority review]